PMDA approval: Viccillin 0.25 g for Injection Viccillin 0.5 g for Injection Viccillin 1 g for Injection Viccillin 2 g for Injection
Applicant: Meiji Seika Pharma Co., Ltd. Approval date: 2012-05-25. Type: Partial change. Active ingredient(s): Ampicillin sodium.
Notes: Drugs with a new additional indication for Listeria monocytogenes as an applicable microorganism and with new additional pediatric and neonate dosages. [Public knowledge-based application after PAFSC's preliminary assessment]